PMDA approval: Arixtra Injection 5 mg Arixtra Injection 7.5 mg
Applicant: GlaxoSmithKline K.K. Approval date: 2011-01-21. Type: New approval. Active ingredient(s): Fondaparinux sodium.
Notes: Drugs with new additional indications and new dosages for the treatment of acute pulmonary thromboembolism and acute deep vein thrombosis.